PMDA approval: Evrysdi Dry Syrup 60 mg
Applicant: Chugai Pharmaceutical Co., Ltd. Approval date: 2024-09-24. Type: Partial change. Active ingredient(s): Risdiplam.
Notes: A drug with a new indication and a new additional pediatric dosage (under 2 months of age) for the treatment of spinal muscular atrophy. [Orphan drug]